PMDA approval: Jemina Tablets
Applicant: Nobelpharma Co., Ltd. Approval date: 2022-03-11. Type: Partial change. Active ingredient(s): Levonorgestrel/ Ethinylestradiol.
Notes: A drug with a new indication and a new dosage for adjusting the start timing of controlled ovarian stimulation in assisted reproductive technology. [Public knowledge-based application]